PMDA approval: Enspryng Syringes for Subcutaneous Injection 120 mg
Applicant: Chugai Pharmaceutical Co., Ltd. Approval date: 2020-06-29. Type: New approval. Active ingredient(s): Satralizumab (genetical recombination).
Notes: A drug with a new active ingredient indicated for the prevention of relapse of neuromyelitis optica spectrum disorder (including neuromyelitis optica). [Orphan drug]